PMDA approval: Rapalimus Tablets 1 mg
Applicant: Nobelpharma Co., Ltd. Approval date: 2014-07-04. Type: New approval. Active ingredient(s): Sirolimus.
Notes: A drug with a new active ingredient indicated for the treatment of lymphangioleiomyomatosis. [Orphan drug]